PMDA approval: Rituxan Intravenous Infusion 100 mg Rituxan Intravenous Infusion 500 mg
Applicant: Zenyaku Kogyo Co., Ltd. Approval date: 2025-03-27. Type: Partial change. Active ingredient(s): Rituximab (genetical recombination).
Notes: Drugs with a new indication and a new dosage for the treatment of nephrotic syndrome (frequently relapsing or steroid-dependent nephrotic syndrome).